PMDA approval: Firazyr Subcutaneous Injection 30 mg Syringe
Applicant: Shire Japan KK. Approval date: 2018-09-21. Type: New approval. Active ingredient(s): Icatibant acetate.
Notes: A drug with a new active ingredient indicated for the treatment of acute attacks of hereditary angioedema. [Orphan drug]